PMDA approval: Vyvdura Combination Subcutaneous Injection
Applicant: Argenx Japan K.K. Approval date: 2024-01-18. Type: New approval. Active ingredient(s): Efgartigimod alfa (genetical recombination) and Vorhyaluronidase alfa (genetical recombination).
Notes: A new combination drug with a new active ingredient indicated for the treatment of generalized myasthenia gravis (for use only in patients who have not sufficiently responded to steroids or other immunosuppressants).